PMDA approval: Enhertu for Intravenous Drip Infusion 100 mg
Applicant: Daiichi Sankyo Company, Limited. Approval date: 2020-09-25. Type: Partial change. Active ingredient(s): Trastuzumab deruxtecan (genetical recombination).
Notes: A drug with a new indication and a new dosage for the treatment of unresectable advanced or recurrent HER2-positive gastric cancer that has progressed after cancer chemotherapy. [SAKIGAKE designation]